PMDA approval: Topotecin Intravenous Drip Infusion 40 mg Topotecin Intravenous Drip Infusion 100 mg
Applicant: Daiichi Sankyo Company, Limited. Approval date: 2013-12-20. Type: Partial change. Active ingredient(s): Irinotecan hydrochloride hydrate.
Notes: Drugs with a new additional indication and a new dosage for the treatment of unresectable pancreatic cancer. [Priority review], [Expedited review]